PMDA approval: Imaavy Intravenous Infusion 300 mg Imaavy Intravenous Infusion 1200 mg
Applicant: Janssen Pharmaceutical K.K. Approval date: 2025-09-19. Type: New approval. Active ingredient(s): Nipocalimab (genetical recombination).
Notes: Drugs with a new active ingredient indicated for the treatment of generalized myasthenia gravis (for use only in patients who have not sufficiently responded to steroids or other immunosuppressants). [Orphan drug]